PMDA approval: Botox for Injection 50 Units Botox for Injection 100 Units
Applicant: GlaxoSmithKline K.K. Approval date: 2019-12-20. Type: Partial change. Active ingredient(s): Botulinum toxin type A.
Notes: Drugs with a new dosage indicated for the treatment of upper limb spasticity.